PMDA approval: Empliciti for I.V. Infusion 300 mg Empliciti for I.V. Infusion 400 mg
Applicant: Bristol-Myers Squibb K.K. Approval date: 2019-11-22. Type: Partial change. Active ingredient(s): Elotuzumab (genetical recombination).
Notes: Drugs with a new dosage indicated for the treatment of relapsed or refractory multiple myeloma. [Orphan drug]